PMDA approval: Abilify Tablets 1 mg Abilify Tablets 3 mg Abilify Tablets 6 mg Abilify Tablets 12 mg Abilify OD Tablets 3 mg Abilify OD Tablets 6 mg Abilify OD Tablets 12 mg Abilify Powder 1% Abilify Oral Solution 0.1%
Applicant: Otsuka Pharmaceutical Co., Ltd. Approval date: 2016-09-28. Type: New approval. Active ingredient(s): Aripiprazole.
Notes: Drugs with a new additional indication, dosage, and dosage form for the treatment of irritability associated with autism spectrum disorder in children and adolescents.